PMDA approval: Lutinus Vaginal Tablet 100 mg
Applicant: Ferring Pharmaceuticals Co.,Ltd. Approval date: 2014-09-26. Type: New approval. Active ingredient(s): Progesterone.
Notes: A drug with a new route of administration indicated for luteal support as part of assisted reproductive technology for infertile women.